PMDA approval: Keytruda Injection 100 mg
Applicant: MSD K.K. Approval date: 2021-11-25. Type: Partial change. Active ingredient(s): Pembrolizumab (genetical recombination).
Notes: A drug with a new indication and a new dosage for the treatment of unresectable advanced or recurrent esophageal cancer.